PMDA approval: Zafatek Tablets 50 mg Zafatek Tablets 100 mg
Applicant: Takeda Pharmaceutical Company Limited. Approval date: 2015-03-26. Type: New approval. Active ingredient(s): Trelagliptin succinate.
Notes: Drugs with a new active ingredient indicated for the treatment of type 2 diabetes mellitus.